PMDA approval: Saphnelo for I.V. Infusion 300 mg
Applicant: AstraZeneca K.K. Approval date: 2021-09-27. Type: New approval. Active ingredient(s): Anifrolumab (genetical recombination).
Notes: A drug with a new active ingredient indicated for the treatment of systemic lupus erythematosus in patients who have not responded sufficiently to conventional treatments.